PMDA approval: Plavix 25 mg Tablets Plavix 75 mg Tablets
Applicant: Sanofi-Aventis K.K. Approval date: 2011-12-22. Type: Partial change. Active ingredient(s): Clopidogrel sulfate.
Notes: Drugs with new additional indications for the treatment of the following ischemic heart disease for which percutaneous coronary intervention (PCI) is applied: stable angina and old myocardial infarction.